PMDA approval: Aimix Combination Tablet LD Aimix Combination Tablet HD
Applicant: Dainippon Sumitomo Pharma Co., Ltd. Approval date: 2012-09-28. Type: New approval. Active ingredient(s): Irbesartan/ amlodipine besilate.
Notes: New combination drugs indicated for the treatment of hypertension.